PMDA approval: Isentress Tablets 600 mg
Applicant: MSD K.K. Approval date: 2018-05-14. Type: New approval. Active ingredient(s): Raltegravir potassium.
Notes: A drug with a new dosage and in an additional dosage form indicated for the treatment of HIV infection. [Orphan drug]